PMDA approval: Venoglobulin IH 5% I.V. 0.5 g/10 mL Venoglobulin IH 5% I.V. 1 g/20 mL Venoglobulin IH 5% I.V. 2.5 g/50 mL Venoglobulin IH 5% I.V. 5 g/100 mL Venoglobulin IH 5% I.V. 10 g/200 mL Venoglobulin IH 10% I.V. 0.5 g/5 mL Venoglobulin IH 10% I.V. 2.5 g/25 mL Venoglobulin IH 10% I.V. 5 g/50 mL Venoglobulin IH 10% I.V. 10 g/100 mL Venoglobulin IH 10% I.V. 20 g/200 mL
Applicant: Japan Blood Products Organization. Approval date: 2019-12-20. Type: Partial change. Active ingredient(s): Polyethylene glycol treated human normal immunoglobulin.
Notes: Drugs with a new additional indication and a new dosage for the preoperative desensitization therapy for kidney transplantation in donor-specific antibodies (DSA) positive patients. [Orphan drug]